PMDA approval: Surepost Tablets 0.25 mg Surepost Tablets 0.5 mg
Applicant: Sumitomo Dainippon Pharma Co., Ltd. Approval date: 2014-11-18. Type: Partial change. Active ingredient(s): Repaglinide.
Notes: Drugs with a revised indication for the treatment of type 2 diabetes mellitus.